PMDA approval: Enosolid Semi Solid for Enteral Use
Applicant: EN Otsuka Pharmaceutical Co., Ltd. Approval date: 2024-09-24. Type: New approval. Active ingredient(s): N/A for this combination drug.
Notes: A combination prescription drug with similar formulations indicated for tube feeding, especially for patients with long-term oral feeding difficulties. It also generally can be used for nutrient retention for postoperative patients.